PMDA approval: Opdivo Intravenous Infusion 240 mg
Applicant: Ono Pharmaceutical Co., Ltd. Approval date: 2018-09-21. Type: New approval. Active ingredient(s): Nivolumab (genetical recombination).
Notes: (1) A drug with new indications and a new dosage in an additional dosage form for the treatment of unresectable or recurrent malignant pleural mesothelioma and melanoma that have progressed after chemotherapy. [Orphan drug] (2) A drug with a new dosage in an additional dosage form indicated for the treatment of melanoma, unresectable or recurrent non-small cell lung cancer, unresectable or metastatic renal cell carcinoma, relapsed or refractory classical Hodgkin lymphoma, recurrent or metastatic head and neck cancer, unresectable or recurrent gastric cancer that has progressed after chemotherapy, and (3) unresectable or metastatic renal cell carcinoma. [(3) Priority review]